PMDA approval: Rituxan Intravenous Infusion 100 mg Rituxan Intravenous Infusion 500 mg
Applicant: Zenyaku Kogyo Co., Ltd. Approval date: 2020-12-25. Type: Partial change. Active ingredient(s): Rituximab (genetical recombination).
Notes: Drugs with a new dosage indicated for the treatment of CD20-positive B-cell non-Hodgkin's lymphoma. [Orphan drug]